PMDA approval: Revolade Tablets 12.5 mg Revolade Tablets 25 mg
Applicant: Novartis Pharma K.K. Approval date: 2017-08-25. Type: Partial change. Active ingredient(s): Eltrombopag olamine.
Notes: Drugs with a new additional indication and a new dosage for the treatment of aplastic anemia. [Orphan drug]